PMDA approval: (1) Infliximab BS for I.V. Infusion 100 mg “NK” (Nippon Kayaku Co., Ltd.) (2) Infliximab BS for I.V. Infusion 100 mg “CTH”
Applicant: Celltrion Inc. Approval date: 2014-07-04. Type: New approval. Active ingredient(s): Infliximab (genetical recombination) [infliximab biosimilar 1].
Notes: Follow-on biologics indicated for the treatment of rheumatoid arthritis in patients who have not responded sufficiently to conventional treatments (including prevention of structural joint damage), the treatment and maintenance therapy of Crohn's disease (for use only in patients in an active phase of moderate to severe Crohn's disease or with external fistula who have not responded sufficiently to conventional treatments), or the treatment of moderate to severe ulcerative colitis (for use only in patients who have not responded sufficiently to conventional treatments).